PMDA approval: Ycanth Topical Solution 0.71%
Applicant: Torii Pharmaceutical Co., Ltd. Approval date: 2025-09-19. Type: New approval. Active ingredient(s): Cantharidin.
Notes: A drug with a new active ingredient indicated for the treatment of molluscum contagiosum.